PMDA approval: Rebetol Capsules 200 mg
Applicant: MSD K.K. Approval date: 2017-03-24. Type: Partial change. Active ingredient(s): Ribavirin.
Notes: A drug with a new additional indication for the improvement of viraemia in patients with chronic hepatitis C or compensated cirrhosis type C in neither Serogroup 1 (genotype 1) nor Serogroup 2 (genotype 2). [Expedited review]